PMDA approval: Darzalex Intravenous Infusion 100 mg Darzalex Intravenous Infusion 400 mg
Applicant: Janssen Pharmaceutical K.K. Approval date: 2020-11-27. Type: Partial change. Active ingredient(s): Daratumumab (genetical recombination).
Notes: Drugs with a new dosage indicated for the treatment of multiple myeloma. [Orphan drug]